PMDA approval: Cerdelga Capsule 100 mg
Applicant: Genzyme Japan K.K. Approval date: 2015-03-26. Type: New approval. Active ingredient(s): Eliglustat tartrate.
Notes: A drug with a new active ingredient indicated for the improvement of various symptoms of Gaucher disease (anemia, thrombocytopenia, hepatosplenomegaly, and bone disease). [Orphan drug]